PMDA approval: Flutiform 50 Aerosol 56 puffs Flutiform 125 Aerosol 56 puffs Flutiform 50 Aerosol 120 puffs Flutiform 125 Aerosol 120 puffs
Applicant: Kyorin Pharmaceutical Co., Ltd. Approval date: 2013-09-20. Type: New approval. Active ingredient(s): (1) Fluticasone propionate (2) Formoterol fumarate hydrate.
Notes: New combination drugs indicated for the treatment of bronchial asthma (when a combination treatment of an inhaled steroid and a long-acting beta-2 agonist is needed).